PMDA approval: Keytruda Injection 20 mg Keytruda Injection 100 mg
Applicant: MSD K.K. Approval date: 2017-11-30. Type: Partial change. Active ingredient(s): Pembrolizumab (genetical recombination).
Notes: Drugs with a new additional indication for the treatment of relapsed or refractory classical Hodgkin's lymphoma.